PMDA approval: Randa Injection 10 mg/20 mL Randa Injection 25 mg/50 mL Randa Injection 50 mg/100 mL
Applicant: Nippon Kayaku Co., Ltd. Approval date: 2024-12-27. Type: Partial change. Active ingredient(s): Cisplatin.
Notes: Drugs with a new indication and a new dosage for the treatment of urothelial carcinoma.